PMDA approval: Noberbar 250 mg for Injection
Applicant: Nobelpharma Co., Ltd. Approval date: 2008-10-16. Type: New approval. Active ingredient(s): Phenobarbital sodium.
Notes: A drug with a new route of administration indicated for the treatment of 1) neonatal seizures and 2) status epilepticus. [Orphan drug: 1]